PMDA approval: Prostandin 20 for Injection
Applicant: Ono Pharmaceutical Co., Ltd. Approval date: 2011-02-23. Type: Partial change. Active ingredient(s): Alprostadil alfadex.
Notes: A drug with a new route of administration and a new dosage for a new additional indication for the diagnosis of erectile disturbance.